PMDA approval: Lumicef Subcutaneous Injection 210 mg Syringe
Applicant: Kyowa Kirin Co., Ltd. Approval date: 2023-08-23. Type: Partial change. Active ingredient(s): Brodalumab (genetical recombination).
Notes: A drug with a new indication for the treatment of palmoplantar pustulosis in patients who have not responded sufficiently to conventional therapies.